PMDA approval: Nesp Injection 5 μg Plastic Syringe Nesp Injection 10 μg Plastic Syringe Nesp Injection 15 μg Plastic Syringe Nesp Injection 20 μg Plastic Syringe Nesp Injection 30 μg Plastic Syringe Nesp Injection 40 μg Plastic Syringe Nesp Injection 60 μg Plastic Syringe Nesp Injection 120 μg Plastic Syringe Nesp Injection 180 μg Plastic Syringe
Applicant: Kyowa Hakko Kirin Co., Ltd. Approval date: 2014-12-18. Type: Partial change. Active ingredient(s): Darbepoetin alfa (genetical recombination).
Notes: Drugs with a new additional indication and a new dosage for the treatment of anemia due to myelodysplastic syndrome. [Orphan drug]